PMDA approval: MyoMIBG-I123 Injection
Applicant: PDRadiopharma Inc. Approval date: 2023-12-22. Type: Partial change. Active ingredient(s): 3-Iodobenzylguanidine (123I).
Notes: A drug with a new indication and a new dosage for cardiac scintigraphy in the diagnoses of Parkinson's disease and dementia with Lewy bodies.